PMDA approval: Evrenzo Tablets 20 mg Evrenzo Tablets 50 mg Evrenzo Tablets 100 mg
Applicant: Astellas Pharma Inc. Approval date: 2019-09-20. Type: New approval. Active ingredient(s): Roxadustat.
Notes: Drugs with a new active ingredient indicated for the treatment of renal anaemia in patients who are undergoing dialysis.